PMDA approval: Vidaza for Injection 100 mg
Applicant: Nippon Shinyaku Co., Ltd. Approval date: 2011-01-21. Type: New approval. Active ingredient(s): Azacitidine.
Notes: A drug with a new active ingredient indicated for the treatment of myelodysplastic syndrome. [Orphan drug]